PMDA approval: Inulead Injection
Applicant: Fuji yakuhin Co., Ltd. Approval date: 2021-08-25. Type: Partial change. Active ingredient(s): Inulin.
Notes: A drug with a new dosage indicated for renal function tests based on measurement of glomerular filtration rate.